PMDA approval: Jardiance Tablets 10 mg
Applicant: Nippon Boehringer Ingelheim Co., Ltd. Approval date: 2024-02-09. Type: Partial change. Active ingredient(s): Empagliflozin.
Notes: A drug with a new indication and a new dosage for the treatment of chronic kidney disease.